PMDA approval: Cymbalta Capsules 20 mg Cymbalta Capsules 30 mg
Applicant: Shionogi & Co., Ltd. Approval date: 2016-03-18. Type: Partial change. Active ingredient(s): Duloxetine hydrochloride.
Notes: Drugs with a new additional indication for the treatment of pain associated with chronic low back pain.